PMDA approval: Nexavar Tablets 200 mg
Applicant: Bayer Yakuhin Ltd. Approval date: 2008-01-25. Type: New approval. Active ingredient(s): Sorafenib tosilate.
Notes: A drug containing a new active ingredient indicated for the treatment of unresectable or metastatic renal cell carcinoma. [Priority review] 1. Zevalin Yttrium (90Y) Injection Set 2. Zevalin Indium (111In) Injection Set (Bayer Yakuhin Ltd.) Approval Approval 1: Ibritumomab tiuxetan (genetical recombination) Yttrium chloride (90Y) 2: Ibritumomab tiuxetan (genetical recombination) Indium chloride (111In) 1: A drug containing a new active ingredient indicated for the treatment of relapsed or refractory, CD20-positive lyphomas of the following: low-grade B-cell non-Hodgkin’s lymphoma and mantle cell lymphoma. 2: A drug containing a new active ingredient indicated for determining the site of ibritumomab tiuxetan (genetical recombination) accumulation. [Orphan drug] Rituxan Injection 10 mg/mL (Zenyaku Kogyo K.K.) Change Rituximab (genetical recombination) Pretreatment for indium (111In) ibritumomab tiuxetan (genetical recombination) injection and yttrium (90Y) ibritumomab tiuxetan (genetical recombination) injection. Expedited review.